PMDA approval: Darzalex Intravenous Infusion 100 mg Darzalex Intravenous Infusion 400 mg
Applicant: Janssen Pharmaceutical K.K. Approval date: 2019-08-22. Type: Partial change. Active ingredient(s): Daratumumab (genetical recombination).
Notes: Drugs with a new indication and a new dosage for the treatment of multiple myeloma. [Orphan drug]